PMDA approval: Tasigna Capsules 150 mg Tasigna Capsules 200 mg
Applicant: Novartis Pharma K.K. Approval date: 2010-12-21. Type: New approval. Active ingredient(s): Nilotinib hydrochloride hydrate.
Notes: Drugs with new additional indications and a new dosage for the treatment of chronic- or accelerated-phase chronic myelocytic leukaemia.